PMDA approval: Insulin Lispro BS Injection HU Solostar "Sanofi" Insulin Lispro BS Injection HU Cart "Sanofi" Insulin Lispro BS Injection HU 100 U/mL "Sanofi"
Applicant: Sanofi K.K. Approval date: 2020-03-25. Type: New approval. Active ingredient(s): Insulin lispro (genetical recombination) [Insulin lispro biosimilar 1].
Notes: Follow-on biologics indicated for the treatment of diabetes mellitus in cases where insulin therapy is indicated.